PMDA approval: Prizbind Intravenous Solution 2.5 g
Applicant: Nippon Boehringer Ingelheim Co., Ltd. Approval date: 2016-09-28. Type: New approval. Active ingredient(s): Idarucizumab (genetical recombination).
Notes: A drug with a new active ingredient indicated for the reversal of anticoagulant effect of dabigatran under the following situations: • In life-threatening or uncontrolled bleeding • When performing emergency surgery or procedures in which serious bleeding is expected. [Priority review]